PMDA approval: Travatanz Ophthalmic Solution 0.004%
Applicant: Alcon Japan Ltd. Approval date: 2007-07-31. Type: New approval. Active ingredient(s): Travoprost.
Notes: A drug containing a new active ingredient indicated for the treatment of glaucoma and ocular hypertension.